PMDA approval: Revcovi 2.4 mg for Intramuscular Injection
Applicant: Teijin Pharma Limited. Approval date: 2019-03-26. Type: New approval. Active ingredient(s): Elapegademase (genetical recombination).
Notes: A drug with a new active ingredient indicated for the treatment of adenosine deaminase deficiency. [Orphan drug]